PMDA approval: Anti-D Globulin for I.M. Injection 1000 "Nichiyaku"
Applicant: Nihon Pharmaceutical Co., Ltd. Approval date: 2011-05-20. Type: Partial change. Active ingredient(s): Dry anti-Rho(D) immune human globulin.
Notes: A drug with a new indication and a new dosage for the prevention of sensitization to the D(Rho) factor in women who are D(Rho) negative and are not already sensitized to the D(Rho) factor, to be used at around 28 weeks of pregnancy or in the case of possible sensitization such as after delivery, miscarriage, artificial abortion, ectopic pregnancy, testing/procedure during pregnancy (e.g., amniocentesis, external cephalic version) or abdominal bruise. [Public knowledge-based application after PAFSC's preliminary assessment]